PMDA approval: Spiramycin Tablets 1.5M IU "Sanofi"
Applicant: Sanofi K.K. Approval date: 2018-07-02. Type: New approval. Active ingredient(s): Spiramycin.
Notes: A drug with a new active ingredient indicated for the prophylaxis of congenital toxoplasmosis. [Orphan drug]